PMDA approval: Actemra 80 mg for Intravenous Infusion Actemra 200 mg for Intravenous Infusion Actemra 400 mg for Intravenous Infusion
Applicant: Chugai Pharmaceutical Co., Ltd. Approval date: 2023-09-25. Type: Partial change. Active ingredient(s): Tocilizumab (genetical recombination).
Notes: Drugs with a new indication for the treatment of cytokine release syndrome induced by the treatment for malignant tumor.